PMDA approval: Darbepoetin Alfa BS Injection 5 μg Syringe "MYL" Darbepoetin Alfa BS Injection 10 μg Syringe "MYL" Darbepoetin Alfa BS Injection 15 μg Syringe "MYL" Darbepoetin Alfa BS Injection 20 μg Syringe "MYL" Darbepoetin Alfa BS Injection 30 μg Syringe "MYL" Darbepoetin Alfa BS Injection 40 μg Syringe "MYL" Darbepoetin Alfa BS Injection 60 μg Syringe "MYL" Darbepoetin Alfa BS Injection 120 μg Syringe "MYL" Darbepoetin Alfa BS Injection 180 μg Syringe "MYL"
Applicant: Mylan EPD G.K. Approval date: 2019-09-20. Type: New approval. Active ingredient(s): Darbepoetin alfa (genetical recombination) [darbepoetin alfa biosimilar 3].
Notes: Follow-on biologics indicated for the treatment of nephrogenic anaemia.